PMDA approval: Ventavis inhalation solution 10 μg
Applicant: Bayer Yakuhin, Ltd. Approval date: 2015-09-28. Type: New approval. Active ingredient(s): Iloprost.
Notes: A drug with a new active ingredient indicated for the treatment of pulmonary arterial hypertension.